PMDA approval: MaQaid Intravitreal Injection 40 mg
Applicant: Wakamoto Pharmaceutical Co., Ltd. Approval date: 2012-11-21. Type: Partial change. Active ingredient(s): Triamcinolone acetonide.
Notes: A drug with a new additional indication and a new dosage for the treatment of diabetic macular edema.